PMDA approval: Racol-NF Semisolid for Enteral Use
Applicant: EN Otsuka Pharmaceutical Co., Ltd. Approval date: 2014-03-24. Type: New approval. Active ingredient(s): N/A for this combination drug.
Notes: A combination prescription drug with similar formulations indicated for tube feeding for especially patients with long-term oral feeding difficulties. It also generally can be used for nutrient retention for postoperative patients.